PMDA approval: Cablivi Injection 10 mg
Applicant: Sanofi K.K. Approval date: 2022-09-26. Type: New approval. Active ingredient(s): Caplacizumab (genetical recombination).
Notes: A drug with a new active ingredient indicated for the treatment of acquired thrombotic thrombocytopenic purpura. [Orphan drug]